PMDA approval: Fosrenol Chewable Tablets 250 mg Fosrenol Chewable Tablets 500 mg Fosrenol Granules 250 mg Fosrenol Granules 500 mg
Applicant: Bayer Yakuhin, Ltd. Approval date: 2013-08-20. Type: Partial change. Active ingredient(s): Lanthanum carbonate hydrate.
Notes: Drugs with a new indication to extend the indication for the improvement of hyperphosphatemia in patients with chronic kidney disease.